PMDA approval: Spikevax Intramuscular Injection
Applicant: Moderna Japan Co., Ltd. Approval date: 2022-09-12. Type: Partial change. Active ingredient(s): Elasomeran Elasomeran and Imelasomeran.
Notes: A drug with a new indication, a new dosage, and other characteristics for the prevention of disease caused by SARS-CoV-2 infection (COVID-19). [Drug pertaining to Special Approval for Emergency]